PMDA approval: Humira 40mg for S.C. Injection Syringe 0.4 mL Humira 40mg for S.C. Injection in pre-filled pen 0.4 mL
Applicant: AbbVie GK. Approval date: 2025-02-20. Type: Partial change. Active ingredient(s): Adalimumab (genetical recombination).
Notes: Drugs with a new indication and a new dosage for the treatment of non-radiographic axial spondyloarthritis in patients who have not responded sufficiently to conventional treatments. [Public knowledge-based application after PAFSC's preliminary assessment]